PMDA approval: Famvir Tab. 250 mg Famciclovir
Applicant: Asahi Kasei Pharma Corporation. Approval date: 2008-04-16. Type: New approval. Active ingredient(s): Famciclovir.
Notes: A drug containing a new active ingredient indicated for the treatment of herpes zoster.